PMDA approval: Datroway for Intravenous Drip Infusion 100 mg
Applicant: Daiichi Sankyo Company, Limited. Approval date: 2024-12-27. Type: New approval. Active ingredient(s): Datopotamab deruxtecan (genetical recombination).
Notes: A drug with a new active ingredient indicated for the treatment of hormone receptor-positive and human epidermal growth factor receptor 2 (HER2)-negative unresectable or recurrent breast cancer in patients who have previously been treated with chemotherapy.